PMDA approval: Taxotere Injection
Applicant: Sanofi-Aventis K.K. Approval date: 2008-08-29. Type: Partial change. Active ingredient(s): Docetaxel hydrate.
Notes: A drug with a new indication and a new dosage for the treatment of prostate cancer. [Priority review]